PMDA approval: Yondelis I.V. infusion 0.25 mg Yondelis I.V. infusion 1 mg
Applicant: Taiho Pharmaceutical Co., Ltd. Approval date: 2015-09-28. Type: New approval. Active ingredient(s): Trabectedin.
Notes: Drugs with a new active ingredient indicated for the treatment of patients with soft tissue sarcoma. [Orphan drug]